PMDA approval: Imjudo Injection 25 mg Imjudo Injection 300 mg
Applicant: AstraZeneca K.K. Approval date: 2022-12-23. Type: New approval. Active ingredient(s): Tremelimumab (genetical recombination).
Notes: Drugs with a new active ingredient indicated for the treatment of unresectable advanced or recurrent non-small-cell lung cancer and unresectable hepatocellular carcinoma.